PMDA approval: Beyfortus 50 mg Solution for Intramuscular Injection in Syringe Beyfortus 100 mg Solution for Intramuscular Injection in Syringe
Applicant: AstraZeneca K.K. Approval date: 2024-03-26. Type: New approval. Active ingredient(s): Nirsevimab (genetical recombination).
Notes: Drugs with a new active ingredient indicated for as follows: 1.The prevention of lower respiratory tract disease caused by respiratory syncytial (RS) virus infection in neonates, infants and children at risk of serious RS virus infection entering their first or second RS virus season after birth. 2.The prophylaxis of lower respiratory tract disease caused by RS virus infection in all neonates and infants other than those described in Item 1 entering their first RS virus season after birth.